PMDA approval: Zyvox Tablets 600 mg Zyvox Injection 600 mg
Applicant: Pfizer Japan Inc. Approval date: 2012-11-21. Type: Partial change. Active ingredient(s): Linezolid.
Notes: Drugs with a new additional pediatric dosage. These drugs are indicated for sepsis, deep skin infection, chronic pyoderma, secondary infection of trauma, burn, and surgical wounds, and pneumonia. [Public knowledge-based application after PAFSC's preliminary assessment]